PMDA approval: Eylea Intravitreal Injection 40 mg/mL Eylea Intravitreal Injection Kit 40 mg/mL
Applicant: Bayer Yakuhin, Ltd. Approval date: 2014-09-19. Type: Partial change. Active ingredient(s): Aflibercept (genetical recombination).
Notes: Drugs with a new additional indication for the treatment of choroidal neovascularization in patients with pathologic myopia.